PMDA approval: Influenza HA Vaccine "Seiken" Flu-Syringe “Seiken”
Applicant: Denka Seiken Co., Ltd. Approval date: 2011-08-08. Type: Partial change. Active ingredient(s): Influenza HA Vaccine.
Notes: Drugs with a new pediatric dosage indicated for the prevention of influenza. [Expedited review]